PMDA approval: 1) Stelara Intravenous Infusion 130 mg 2) Stelara Subcutaneous Injection 45 mg Syringe
Applicant: Janssen Pharmaceutical K.K. Approval date: 2020-03-25. Type: Partial change. Active ingredient(s): Ustekinumab (genetical recombination).
Notes: 1) A drug with a new indication for the remission induction therapy of moderate to severe ulcerative colitis (for use only in patients who have not sufficiently responded to conventional treatments). 2) A drug with a new indication for the maintenance therapy of moderate to severe ulcerative colitis (for use only in patients who have not sufficiently responded to conventional treatments).